PMDA approval: Prograf Capsules 0.5 mg Prograf Capsules 1 mg Prograf Capsules 5 mg Prograf Granules 0.2 mg Prograf Granules 1 mg Graceptor Capsules 0.5 mg Graceptor Capsules 1 mg Graceptor Capsules 5 mg
Applicant: Astellas Pharma Inc. Approval date: 2023-12-22. Type: Partial change. Active ingredient(s): Tacrolimus hydrate.
Notes: Drugs with a new dosage indicated for the suppression of rejection reaction associated with kidney transplantation. [Orphan drug]